PMDA approval: Vyloy for I.V. Infusion 100 mg
Applicant: Astellas Pharma Inc. Approval date: 2024-03-26. Type: New approval. Active ingredient(s): Zolbetuximab (genetical recombination).
Notes: A drug with a new active ingredient indicated for the treatment of CLDN18.2 (Claudin-18 splice variant 2)-positive unresectable advanced or recurrent gastric cancer.